PMDA approval: Oblean Tablets 120 mg
Applicant: Takeda Pharmaceutical Company Limited. Approval date: 2013-09-20. Type: New approval. Active ingredient(s): Cetilistat.
Notes: A drug with a new active ingredient indicated for the treatment of obesity (for use only in patients who have both type 2 diabetes mellitus and dyslipidaemia and whose BMI level is 25kg/m 2 or more even with diet and exercise therapies).